PMDA approval: Toviaz Tablet 4 mg Toviaz Tablet 8mg
Applicant: Pfizer Japan Inc. Approval date: 2012-12-25. Type: New approval. Active ingredient(s): Fesoterodine fumarate.
Notes: Drugs with a new active ingredient indicated for the treatment of urinary urgency, urinary frequency, and urge urinary incontinence associated with overactive bladder. 62 (1) Takepron capsules 15 Takepron capsules 30 (2) Takepron OD Tablets 15 Takepron OD Tablets 30 (Takeda Pharmaceutical Company Limited) (3) Omepral Tablets 10 Omepral Tablets 20 (AstraZeneca K.K.) (4) Omeprazon Tablets 10 mg Omeprazon Tablets 20 mg (Mitsubishi Tanabe Pharma Corporation) (5) Omeprazole Tablets 10 mg "Mylan" Omeprazole Tablets 20 mg "Mylan" (Mylan Seiyaku Ltd.) (6) Pariet Tablets 10 mg (Eisai Co., Ltd.) (7) Rabeprazole Na Tablets 10 mg "Ohara" (Ohara Pharmaceutical Co., Ltd.) (8) Nexium Capsules 10 mg Nexium Capsules 20 mg (AstraZeneca K.K.) Change Change Change Change Change Change Change Change Change Change Change Change Change Change (1)(2) Lansoprazole (3)(4)(5) Omeprazole (6)(7) Rabeprazole sodium (8) Esomeprazole magnesium hydrate (9)(10)(11)(12)(13)(14) Amoxicillin hydrate (15)(16)(17)(18) Clarithromycin (19) Metronidazole (20) Lansoprazole/amoxicillin hydrate/clarithromycin